PMDA approval: Ultibro Inhalation Capsules
Applicant: Novartis Pharma K.K. Approval date: 2013-09-20. Type: New approval. Active ingredient(s): (1) Indacaterol maleate (2) Glycopyrronium bromide.
Notes: A new combination drug indicated for the relief of symptoms secondary to airway obstructive disorder in chronic obstructive pulmonary disease (chronic bronchitis, emphysema) (when a combination treatment of an inhaled long-acting anticholinergic and a long-acting beta-2 agonist is needed).